PMDA approval: Gemzar Injection 200 mg Gemzar Injection 1 g (Eli Lilly Japan K.K.) Gemcitabine for I.V. infusion 200 mg "Yakult" Gemcitabine for I.V. infusion 1 g "Yakult" (Takata Seiyaku Co., Ltd.) Gemcitabine for i.v. infusion 200 mg "Sawai" Gemcitabine for i.v. infusion 1 g "Sawai" (Sawai Pharmaceutical Co., Ltd.) Gemcitabine for I.V. Infusion 200 mg "NK" Gemcitabine for I.V. Infusion 1 g "NK" (Nippon Kayaku Co., Ltd.) Gemcitabine for I.V. Infusion 200 mg "Hospira" Gemcitabine for I.V. Infusion 1 g "Hospira"
Applicant: Hospira Japan Co., Ltd. Approval date: 2013-02-21. Type: Partial change. Active ingredient(s): Gemcitabine hydrochloride.
Notes: Drugs with a new additional indication and a new dosage for the treatment of relapsed or refractory malignant lymphoma. [Public knowledge-based application after PAFSC's preliminary assessment]